PMDA approval: Verzenio Tablets 50 mg Verzenio Tablets 100 mg Verzenio Tablets 150 mg
Applicant: Eli Lilly Japan K.K. Approval date: 2021-12-24. Type: Partial change. Active ingredient(s): Abemaciclib.
Notes: Drugs with a new indication and a new dosage for the postoperative adjuvant treatment of hormone receptor (HR)-positive and human epidermal growth factor receptor 2 (HER2)-negative breast cancer with a high risk of recurrence.